PMDA approval: E Keppra Dry Syrup 50% E Keppra for I.V. Infusion 500 mg
Applicant: UCB Japan Co., Ltd. Approval date: 2023-06-26. Type: Partial change. Active ingredient(s): Levetiracetam.
Notes: Drugs with a new additional pediatric dosage indicated for the treatment of partial seizures (including secondary generalized seizures) in patients with epilepsy.